PMDA approval: Ovidrel Syringe 250 μg
Applicant: Merck Serono Co., Ltd. Approval date: 2016-09-28. Type: New approval. Active ingredient(s): Choriogonadotropin alfa (genetical recombination).
Notes: A drug with a new active ingredient indicated for: • Induction of ovulation and luteinization in patients with anovulation or oligoovulation associated with hypothalamic-pituitary dysfunction • Induction of final follicular maturation and luteinization as part of assisted reproductive technology for infertile women.